PMDA approval: Edurant Tablets 25 mg
Applicant: Janssen Pharmaceutical K.K. Approval date: 2012-05-18. Type: New approval. Active ingredient(s): Rilpivirine hydrochloride.
Notes: A drug with a new active ingredient indicated for the treatment of HIV-1 infection. [Orphan drug]